PMDA approval: Sutent Capsule 12.5 mg
Applicant: Pfizer Japan Inc. Approval date: 2008-04-16. Type: New approval. Active ingredient(s): Sunitinib malate.
Notes: A drug containing a new active ingredient indicated for the treatment of imatinib-resistant gastrointestinal stromal tumors, and unresectable or metastatic renal cell carcinomas. [Priority review]